PMDA approval: Onoact for Intravenous Infusion 50 mg Onoact for Intravenous Infusion 150 mg
Applicant: Ono Pharmaceutical Co., Ltd. Approval date: 2020-06-29. Type: Partial change. Active ingredient(s): Landiolol hydrochloride.
Notes: Drugs with a new indication and a new dosage for the treatment of the following tachyarrhythmia associated with sepsis: atrial fibrillation, atrial flutter, sinus tachycardia.